PMDA approval: Alesion Ophthalmic Solutoin 0.05%
Applicant: Santen Pharmaceutical Co., Ltd. Approval date: 2013-09-20. Type: New approval. Active ingredient(s): Epinastine hydrochloride.
Notes: A drug with a new route of administration indicated for the treatment of allergic conjunctivitis.